PMDA approval: Squarekids Subcutaneous Injection Syringe
Applicant: Kitasato Daiichi Sankyo Vaccine Co., Ltd. Approval date: 2014-07-04. Type: New approval. Active ingredient(s): Adsorbed diphtheria-purified pertussis-tetanus-inactivated polio (salk vaccine) combined vaccine.
Notes: A new combination drug indicated for the prevention of pertussis, diphtheria, tetanus, and acute poliomyelitis.